PMDA approval: Musredo Tablets 5 mg Musredo Tablets 12.5 mg Musredo Tablets 25 mg Musredo Tablets 50 mg Musredo Tablets 75 mg
Applicant: Bayer Yakuhin, Ltd. Approval date: 2021-01-22. Type: New approval. Active ingredient(s): Molidustat sodium.
Notes: Drugs with a new active ingredient indicated for the treatment of renal anemia.